PMDA approval: Selara Tablets 25 mg Selara Tablets 50 mg
Applicant: Pfizer Japan Inc. Approval date: 2016-12-19. Type: Partial change. Active ingredient(s): Eplerenone.
Notes: Drugs with a new additional indication and a new dosage for the treatment of chronic heart failure in patients receiving basic treatment with angiotensin converting enzyme inhibitors or angiotensin II receptor antagonists, β-blockers, diuretics, etc.